PMDA approval: Alecensa Capsule 20 mg Alecensa Capsule 40 mg
Applicant: Chugai Pharmaceutical Co., Ltd. Approval date: 2014-07-04. Type: New approval. Active ingredient(s): Alectinib hydrochloride.
Notes: Drugs with a new active ingredient indicated for the treatment of unresectable advanced/relapsed ALK fusion gene-positive non-small-cell lung cancer. [Orphan drug]